PMDA approval: (1) Skyrizi Intravenous Infusion 600 mg (2) Skyrizi for Subcutaneous Injection 360 mg Auto Dosers
Applicant: AbbVie GK. Approval date: 2022-09-26. Type: New approval. Active ingredient(s): Risankizumab (genetical recombination).
Notes: (1) A drug with a new route of administration indicated for the remission induction therapy of moderate to severe active Crohn’s disease (for use only in patients who have not sufficiently responded to conventional treatments). (2) A drug with a new indication and a new dosage in an additional dosage form for the maintenance therapy of moderate to severe active Crohn’s disease (for use only in patients who have not sufficiently responded to conventional treatments).